PMDA approval: Malarone Combination Tablets
Applicant: GlaxoSmithKline K.K. Approval date: 2012-12-25. Type: New approval. Active ingredient(s): Atovaquone/proguanil hydrochloride.
Notes: A new combination drug with a new active ingredient indicated for the treatment and prevention of malaria. [Priority review]